PMDA approval: Poteligeo Injection 20 mg
Applicant: Kyowa Hakko Kirin Co., Ltd. Approval date: 2018-08-21. Type: Partial change. Active ingredient(s): Mogamulizumab (genetical recombination).
Notes: A drug with new indications and a new dosage for the treatment of relapsed or refractory cutaneous T-cell lymphoma. [Orphan drug]